PMDA approval: Duvroq Tablets 1 mg Duvroq Tablets 2 mg Duvroq Tablets 4 mg Duvroq Tablets 6 mg
Applicant: GlaxoSmithKline K.K. Approval date: 2020-06-29. Type: New approval. Active ingredient(s): Daprodustat.
Notes: Drugs with a new active ingredient indicated for the treatment of renal anemia.